PMDA approval: Calonal Tab. 500
Applicant: Showa Yakuhin Kako Co., Ltd. Approval date: 2014-09-26. Type: New approval. Active ingredient(s): Acetaminophen.
Notes: A drug in an additional dosage form. Note: the product was submitted for an approval application as a drug with a new additional indication for the treatment of osteoarthrosis and with an expanded dosage of acetaminophen in an additional dosage form. The new additional indication and the expanded dosage were approved on January 21, 2011, whereas approval of the additional dosage form had been under review.